PMDA approval: Calquence Capsules 100 mg
Applicant: AstraZeneca K.K. Approval date: 2022-12-23. Type: Partial change. Active ingredient(s): Acalabrutinib.
Notes: A drug with a new indication for the treatment of chronic lymphocytic leukemia (including small lymphocytic lymphoma).